PMDA approval: Byetta Subcutaneous Injection 5 μg Pen 300 Byetta Subcutaneous Injection 10 μg Pen 600 Byetta Subcutaneous Injection 10 μg Pen 300
Applicant: Eli Lilly Japan K.K. Approval date: 2010-10-27. Type: New approval. Active ingredient(s): Exenatide.
Notes: Drugs with a new active ingredient indicated for the treatment of type 2 diabetes mellitus (for use only in patients who have not responded sufficiently to treatment with sulfinylurea [including concomitant treatment with biguanides or thiazolidines] along with diet and exercise therapies).